PMDA approval: Votrient Tablets 200 mg
Applicant: GlaxoSmithKline K.K. Approval date: 2014-03-17. Type: Partial change. Active ingredient(s): Pazopanib hydrochloride.
Notes: A drug with a new additional indication for the treatment of unresectable or metastatic renal cell carcinoma.